PMDA approval: Isovorin Injection 25 mg Isovorin Injection 100 mg
Applicant: Pfizer Japan Inc. Approval date: 2023-03-27. Type: Partial change. Active ingredient(s): Calcium levofolinate hydrate.
Notes: Drugs with a new indication and a new dosage for the treatment of unresectable advanced or recurrent gastric cancer. [Public knowledge-based application after PAESC's preliminary assessment]